PMDA approval: Jevtana 60 mg I.V. Infusion
Applicant: Sanofi K.K. Approval date: 2014-07-04. Type: New approval. Active ingredient(s): Cabazitaxel acetonate.
Notes: A drug with a new active ingredient indicated for the treatment of prostate cancer. [Priority review]